PMDA approval: Cosentyx for S.C. Injection 150 mg Syringe Cosentyx for S.C. Injection 150 mg Pen
Applicant: Novartis Pharma K.K. Approval date: 2020-08-21. Type: Partial change. Active ingredient(s): Secukinumab (genetical recombination).
Notes: Drugs with a new indication and a new dosage for the treatment of non-radiographic axial spondyloarthritis in patients who have not responded sufficiently to conventional treatments. [Orphan drug]